PMDA approval: Beyonttra Tablets 400 mg
Applicant: Alexion Pharma GK. Approval date: 2025-03-27. Type: New approval. Active ingredient(s): Acoramidis hydrochloride.
Notes: A drug with a new active ingredient indicated for the treatment of transthyretin cardiac amyloidosis (wild-type and hereditary).